PMDA approval: Rituxan Intravenous Infusion 100 mg Rituxan Intravenous Infusion 500 mg
Applicant: Zenyaku Kogyo Co., Ltd. Approval date: 2022-06-20. Type: Partial change. Active ingredient(s): Rituximab (genetical recombination).
Notes: Drugs with a new indication and a new dosage for the prevention of relapse of neuromyelitis optica spectrum disorder (including neuromyelitis optica). [Orphan drug]